PMDA approval: Hycamtin for Injection 1.1 mg
Applicant: Nippon Kayaku Co., Ltd. Approval date: 2015-11-20. Type: Partial change. Active ingredient(s): Nogitecan hydrochloride.
Notes: A drug with a new additional indication and a new dosage for the treatment of advanced or recurrent cervical cancer.